PMDA approval: Taxol Injection 30 mg Taxol Injection 100 mg (Bristol-Myers K.K.) Paclitaxel Inj. 30 mg/5 mL 'NK' Paclitaxel Inj. 100 mg/16.7 mL 'NK' (Nippon Kayaku Co., Ltd.) Paclitaxel Injection 30 mg 'Sawai' Paclitaxel Injection 100 mg 'Sawai' Paclitaxel Injection 150 mg 'Sawai' (Sawai Pharmaceutical Co., Ltd.) Paclitaxel Intravenous Infusion 30 mg 'Sandoz' Paclitaxel Intravenous Infusion 100 mg 'Sandoz'
Applicant: Sandoz K.K. Approval date: 2012-03-21. Type: Partial change. Active ingredient(s): Paclitaxel.
Notes: Drugs with new additional indications and new dosages for the treatment of relapsed or metastatic head and neck cancer, relapsed or metastatic esophagus cancer, angiosarcoma, advanced or relapsed cervical cancer. A new dosage for once-weekly administration for ovarian cancer has also been added. [Public knowledge-based application after PAFSC's preliminary assessment]